PMDA approval: Flagyl Oral Tablet 250 mg
Applicant: Shionogi & Co., Ltd. Approval date: 2012-08-10. Type: Partial change. Active ingredient(s): Metronidazole.
Notes: A drug with new additional indications and a new dosage for the treatment of anaerobic bacterial infection, infectious enteritis, amoebic dysentery, and Giardia lamblia infection. [Public knowledge-based application after PAFSC's preliminary assessment]